PMDA approval: Otezla Tablets 10 mg Otezla Tablets 20 mg Otezla Tablets 30 mg
Applicant: Celgene K.K. Approval date: 2019-09-20. Type: Partial change. Active ingredient(s): Apremilast.
Notes: Drugs with a new indication for the treatment of oral ulcer associated with Behcet’s disease in patients who have not responded sufficiently to local treatment.